PMDA approval: Cystadrops Ophthalmic Solution 0.38%
Applicant: Viatris Pharmaceuticals Japan Inc. Approval date: 2024-03-26. Type: New approval. Active ingredient(s): Cysteamine hydrochloride.
Notes: A drug with a new active ingredient indicated for the reduction of corneal cystine crystals in patients with cystinosis. [Orphan drug]